PMDA approval: Rinderon Injection 2 mg (0.4%) Rinderon Injection 4 mg (0.4%)
Applicant: Shionogi & Co., Ltd. Approval date: 2009-11-06. Type: Partial change. Active ingredient(s): Betamethasone sodium phosphate.
Notes: Drugs with a new additional indication and a new dosage for prevention of neonatal respiratory distress syndrome by way of enhancing fetal lung maturation by maternal administration for use in cases where premature birth is expected.